PMDA approval: Vectibix for Intravenous Infusion 100 mg (Takeda Pharmaceutical Company Limited) Vectibix for Intravenous Infusion 100 mg "Takeda Bio"
Applicant: Takeda Bio Development Center Limited. Approval date: 2010-04-16. Type: New approval. Active ingredient(s): Panitumumab (genetical recombination）.
Notes: Drugs with a new active ingredient indicated for the treatment of unresectable, advanced or recurrent colorectal cancer with wild-type KRAS.